PMDA approval: Spydia Nasal Spray 5 mg Spydia Nasal Spray 7.5 mg Spydia Nasal Spray 10 mg
Applicant: Aculys Pharma, Inc. Approval date: 2025-06-24. Type: New approval. Active ingredient(s): Diazepam.
Notes: Drugs with a new route of administration indicated for the treatment of status epilepticus. [Orphan drug]